PMDA approval: Aloxi I.V. Injection 0.75 mg Aloxi I.V. Infusion Bag 0.75 mg
Applicant: Taiho Pharmaceutical Co., Ltd. Approval date: 2021-05-27. Type: Partial change. Active ingredient(s): Palonosetron hydrochloride.
Notes: Drugs with a new additional pediatric dosage indicated for the treatment of digestive symptoms (nausea and vomiting, including delayed phase) resulting from the administration of antineoplastic agents (cisplatin, etc.).